PMDA approval: Kisunla Intravenous Infusion 350 mg
Applicant: Eli Lilly Japan K.K. Approval date: 2024-09-24. Type: New approval. Active ingredient(s): Donanemab (genetical recombination).
Notes: A drug with a new active ingredient indicated for slowing the progression of mild cognitive impairment and mild dementia due to Alzheimer’s disease.